PMDA approval: Zintus Tablets 25 mg Zintus Tablets 50 mg
Applicant: Nobelpharma Co., Ltd. Approval date: 2024-03-26. Type: New approval. Active ingredient(s): Zinc histidine hydrate.
Notes: Drugs with a new active ingredient indicated for the treatment of hypozincemia.